PMDA approval: Duphaston Tablets 5mg
Applicant: Mylan EPD G.K. Approval date: 2022-03-11. Type: Partial change. Active ingredient(s): Dydrogesterone.
Notes: A drug with a new indication and a new dosage for the prevention of premature ovulation during controlled ovarian stimulation. [Public knowledge-based application]